PMDA approval: Revlimid Capsules 2.5 mg Revlimid Capsules 5 mg
Applicant: Bristol-Myers Squibb K.K. Approval date: 2025-02-20. Type: Partial change. Active ingredient(s): Lenalidomide hydrate.
Notes: Drugs with a new dosage indicated for the treatment of multiple myeloma.